PMDA approval: Longes Tablets 5 mg Longes Tablets 10 mg Longes Tablets 20 mg
Applicant: Shionogi & Co., Ltd. Approval date: 2012-06-22. Type: Partial change. Active ingredient(s): Lisinopril hydrate.
Notes: Drugs with a new additional pediatric dosage. These drugs are indicated for the treatment of hypertension. [Public knowledge-based application after PAFSC's preliminary assessment] 2 Jun.22, 2012 17 Norvasc Tablets 2.5 mg Norvasc Tablets 5 mg Norvasc OD Tablets 2.5 mg Norvasc OD Tablets 5 mg (Pfizer Japan Inc.) Amlodin Tablets 2.5 mg Amlodin Tablets 5 mg Amlodin OD Tablets 2.5 mg Amlodin OD Tablets 5 mg (Dainippon Sumitomo Pharma Co., Ltd.) Change Change Change Change Change Change Change Change Amlodipine besilate Drugs with a new additional pediatric dosage. These drugs are indicated for the treatment of hypertension. [Public knowledge-based application after PAFSC's preliminary assessment]